PMDA approval: Radiogardase Capsule 500 mg
Applicant: Nihon Medi-Physics Co., Ltd. Approval date: 2012-12-21. Type: Partial change. Active ingredient(s): Iron (III) hexacyanoferrate (II).
Notes: A drug with a new additional indication for the treatment of thallium and thallium compound poisoning.